PMDA approval: Lagevrio Capsules 200 mg
Applicant: MSD K.K. Approval date: 2021-12-24. Type: New approval. Active ingredient(s): Molnupiravir.
Notes: A drug with a new active ingredient indicated for the treatment of disease caused by SARS-CoV-2 infection (COVID-19). [Drug pertaining to Special Approval for Emergency]